PMDA approval: Ciproxan-I.V. 200
Applicant: Bayer Yakuhin, Ltd. Approval date: 2015-09-24. Type: Partial change. Active ingredient(s): Ciprofloxacin.
Notes: A drug with a new increased dose for adults indicated for the treatment of sepsis, pneumonia, etc., and also with new indications and new dosages for children for both the treatment of complicated cystitis, pyelonephritis and anthrax, and the improvement of symptoms associated with respiratory infection caused by Pseudomonas aeruginosa in cystic fibrosis.